PMDA approval: Camzyos Capsules 5 mg Camzyos Capsules 2.5 mg Camzyos Capsules 1 mg
Applicant: Bristol-Myers Squibb K.K. Approval date: 2025-03-27. Type: New approval. Active ingredient(s): Mavacamten.
Notes: Drugs with a new active ingredient indicated for the treatment of symptomatic obstructive hypertrophic cardiomyopathy. [Orphan drug]